PMDA approval: Vaxem Hib Suspension Liquid for Injection
Applicant: Takeda Pharmaceutical Company Limited. Approval date: 2016-01-22. Type: New approval. Active ingredient(s): Haemophilus influenzae type b vaccine absorbed (mutated diphtheria CRM197 conjugate).
Notes: A drug with a new active ingredient indicated for the prophylaxis of Haemophilus influenzae type b infections.